PMDA approval: Ronapreve for Intravenous Infusion Set 300 Ronapreve for Intravenous Infusion Set 1332
Applicant: Chugai Pharmaceutical Co., Ltd. Approval date: 2021-11-05. Type: Partial change. Active ingredient(s): Casirivimab (genetical recombination), Imdevimab (genetical recombination).
Notes: Drugs with a new route of administration and a new indication for the treatment and suppression of development of disease caused by SARS-CoV- 2 infection (COVID-19). [Drug pertaining to Special Approval for Emergency]